PMDA approval: Rituximab BS Intravenous Infusion 100 mg [Pfizer] Rituximab BS Intravenous Infusion 500 mg [Pfizer]
Applicant: Pfizer Japan Inc. Approval date: 2019-09-20. Type: New approval. Active ingredient(s): Rituximab (genetical recombination) [rituximab biosimilar 2].
Notes: Follow-on biologics indicated for the treatment of CD20-positive B-cell non-Hodgkin’s lymphoma, CD20-positive B-cell lymphoproliferative disorder associated with immunosuppression, granulomatosis with polyangiitis, and microscopic polyangiitis.